PMDA approval: Rinvoq Tablets 30 mg
Applicant: AbbVie GK. Approval date: 2021-09-27. Type: New approval. Active ingredient(s): Upadacitinib hydrate.
Notes: A drug in an additional dosage form indicated for the treatment of atopic dermatitis in patients who have not responded sufficiently to conventional treatments.